PMDA approval: Treakisym Injection 25 mg Treakisym Injection 100 mg
Applicant: SymBio Pharmaceuticals Limited. Approval date: 2018-07-02. Type: Partial change. Active ingredient(s): Bendamustine hydrochloride.
Notes: Drugs with a new dosage indicated for the treatment of low-grade B-cell non-Hodgkin’s lymphoma.